PMDA approval: Trerief Tablets 25 mg Trerief OD Tablets 25 mg
Applicant: Sumitomo Dainippon Pharma Co., Ltd. Approval date: 2018-07-02. Type: Partial change. Active ingredient(s): Zonisamide.
Notes: Drugs with a new additional indication and a new dosage for the treatment of parkinsonism in patients with dementia with Lewy bodies (in the cases where parkinsonism persists after using levodopa-containing products).